PMDA approval: Fintepla Oral Solution 2.2 mg/mL
Applicant: UCB Japan Co., Ltd. Approval date: 2024-03-26. Type: Partial change. Active ingredient(s): Fenfluramine hydrochloride.
Notes: A drug with a new indication and a new dosage for use as an adjunctive therapy with other antiepileptic drugs to treat epileptic seizures in patients with Lennox-Gastaut syndrome who have not responded sufficiently to other antiepileptic drugs. [Orphan drug]